PMDA approval: Radicut Inj. 30 mg Radicut Bag for I.V. Infusion 30 mg
Applicant: Mitsubishi Tanabe Pharma Corporation. Approval date: 2015-06-26. Type: Partial change. Active ingredient(s): Edaravone.
Notes: Drugs with a new additional indication and a new dosage for delaying the functional disorder in patients with amyotrophic lateral sclerosis (ALS). [Orphan drug]